PMDA approval: Lipacreon Granules 300 mg Sachet Lipacreon Capsules 150 mg
Applicant: Abbott Japan Co., Ltd. Approval date: 2011-04-22. Type: New approval. Active ingredient(s): Pancrelipase.
Notes: Drugs with a new active ingredient indicated for the replacement of pancreatic digestive enzymes in pancreatic exocrine insufficiency.